PMDA approval: Imfinzi Injection 120 mg Imfinzi Injection 500 mg
Applicant: AstraZeneca K.K. Approval date: 2022-12-23. Type: Partial change. Active ingredient(s): Durvalumab (genetical recombination).
Notes: (1) Drugs with a new indication and a new dosage for the treatment of unresectable advanced or recurrent non-small-cell lung cancer. (2) Drugs with a new indication and a new dosage for the treatment of unresectable hepatocellular carcinoma.